PMDA approval: Endoxan for Injection 100 mg Endoxan for Injection 500 mg Endoxan Tablets 50 mg
Applicant: Shionogi & Co., Ltd. Approval date: 2011-02-23. Type: Partial change. Active ingredient(s): Cyclophosphamide.
Notes: Drugs with a new additional indication and new dosages for treatment-resistant rheumatic disease in adults and children. [Public knowledge-based application after PAFSC's preliminary assessment]